PMDA approval: Keytruda Injection 100 mg
Applicant: MSD K.K. Approval date: 2022-02-25. Type: Partial change. Active ingredient(s): Pembrolizumab (genetical recombination).
Notes: A drug with a new dosage indicated for the treatment of unresectable or metastatic renal cell carcinoma.